PMDA approval: Certican Tablets 0.25 mg Certican Tablets 0.5 mg Certican Tablets 0.75 mg
Applicant: Novartis Pharma K.K. Approval date: 2011-12-22. Type: Partial change. Active ingredient(s): Everolimus.
Notes: Drugs with a new additional indication and a new dosage for the inhibition of rejection in renal transplantation.